PMDA approval: Victoza Subcutaneous Injection 18 mg
Applicant: Novo Nordisk Pharma Ltd. Approval date: 2019-05-22. Type: Partial change. Active ingredient(s): Liraglutide (genetical recombination).
Notes: A drug with a new dosage and other characteristics indicated for the treatment of type 2 diabetes mellitus.